PMDA approval: Avastin 100 mg/4 mL Intravenous Infusion Avastin 400 mg/16 mL Intravenous Infusion
Applicant: Chugai Pharmaceutical Co., Ltd. Approval date: 2013-06-14. Type: Partial change. Active ingredient(s): Bevacizumab (genetical recombination).
Notes: Drugs with a new additional indication and a new dosage for the treatment of malignant glioma. [Orphan drug]